PMDA approval: Jaypirca Tablets 50 mg Jaypirca Tablets 100 mg
Applicant: Eli Lilly Japan K.K. Approval date: 2024-06-24. Type: New approval. Active ingredient(s): Pirtobrutinib.
Notes: Drugs with a new active ingredient indicated for the treatment of relapsed or refractory mantle cell lymphoma with resistance or intolerance to other BTK inhibitors.